PMDA approval: HyQvia 10% S.C. Injection Set 5 g/50 mL HyQvia 10% S.C. Injection Set 10 g/100 mL HyQvia 10% S.C. Injection Set 20 g/200 mL
Applicant: Takeda Pharmaceutical Company Limited. Approval date: 2024-12-27. Type: New approval. Active ingredient(s): Human normal immunoglobulin G (pH4-treated acidic normal human immunoglobulin [subcutaneous injection]) and Vorhyaluronidase alfa (genetical recombination).
Notes: Drugs with a new active ingredient indicated for the treatment of agammaglobulinemia or hypogammaglobulinemia.